PMDA approval: Afinitor Dispersible Tablet 2 mg Afinitor Dispersible Tablet 3 mg
Applicant: Novartis Pharma K.K. Approval date: 2012-12-25. Type: New approval. Active ingredient(s): Everolimus.
Notes: Drugs with a new additional indication and a new dosage in an additional dosage form for the treatment of subependymal giant cell astrocytoma associated with tuberous sclerosis complex (currently in the reexamination period). [Orphan drug]